PMDA approval: Leqvio for S.C. Injection Syringe 300 mg
Applicant: Novartis Pharma K.K. Approval date: 2023-09-25. Type: New approval. Active ingredient(s): Inclisiran sodium.
Notes: A drug with a new active ingredient indicated for the treatment of familial hypercholesterolemia and hypercholesterolemia (for use only in patients who met all of the followings:) • High risk of developing cardiovascular events • Patients who have not sufficiently responded to an HMG-CoA reductase inhibitor or in whom treatment with HMG-CoA reductase inhibitors is not suitable.